PMDA approval: Kcentra for I.V. Injection 500 Kcentra for I.V. Injection 1000
Applicant: CSL Behring K.K. Approval date: 2017-03-30. Type: New approval. Active ingredient(s): Freeze-dried human prothrombin complex concentrated.
Notes: Drugs with a new active ingredient indicated for the control of bleeding tendency in patients treated with vitamin K antagonists at the time of acute serious bleeding or an emergency surgery/procedures in which serious bleeding is expected. [Orphan drug]